PMDA approval: Suglat Tablets 25 mg Suglat Tablets 50 mg
Applicant: Astellas Pharma Inc. Approval date: 2018-12-21. Type: Partial change. Active ingredient(s): Ipragliflozin L-proline.
Notes: Drugs with a new additional indication and a new dosage for the treatment of type 1 diabetes mellitus.